PMDA approval: Opdivo Intravenous Infusion 20 mg Opdivo Intravenous Infusion 100 mg
Applicant: Ono Pharmaceutical Co., Ltd. Approval date: 2014-07-04. Type: New approval. Active ingredient(s): Nivolumab (genetical recombination).
Notes: Drugs with a new active ingredient indicated for the treatment of unresectable malignant melanoma. [Orphan drug]